PMDA approval: Yaz Flex Combination Tablets
Applicant: Bayer Yakuhin, Ltd. Approval date: 2022-03-11. Type: Partial change. Active ingredient(s): Drospirenone/ Ethinylestradiol betadex.
Notes: A drug with a new indication and a new dosage for adjusting the start timing of controlled ovarian stimulation in assisted reproductive technology. [Public knowledge-based application]